PMDA approval: Velsipity Tablets 2 mg
Applicant: Pfizer Japan Inc. Approval date: 2025-06-24. Type: New approval. Active ingredient(s): Etrasimod L-arginine.
Notes: A drug with a new active ingredient indicated for the treatment of moderate to severe ulcerative colitis (for use only in patients who have not sufficiently responded to conventional treatments).